PMDA approval: Diagnogreen for Injection 25 mg
Applicant: Daiichi Sankyo Company, Limited. Approval date: 2012-02-22. Type: Partial change. Active ingredient(s): Indocyanine green.
Notes: A drug with a new additional indication and a new dosage for the use of cerebral angiography (by fluorescence measurement in infrared-ray irradiation) in neurosurgical operation. [Public knowledge-based application after PAFSC's preliminary assessment]